PMDA approval: Hysron Tablets 5
Applicant: Kyowa Kirin Co., Ltd. Approval date: 2022-03-11. Type: Partial change. Active ingredient(s): Medroxyprogesterone acetate.
Notes: A drug with a new indication and a new dosage for the prevention of premature ovulation during controlled ovarian stimulation. [Public knowledge-based application]